PMDA approval: Rituxan Injection 10 mg/mL
Applicant: Zenyaku Kogyo Co., Ltd. Approval date: 2014-08-29. Type: Partial change. Active ingredient(s): Rituximab (genetical recombination).
Notes: A drug with a new additional indication and a new dosage for the treatment of refractory nephrotic syndrome (for use in patients with frequent recurrence or steroid-dependent). [Orphan drug]